PMDA approval: Abraxane I.V. Infusion 100 mg
Applicant: Taiho Pharmaceutical Co., Ltd. Approval date: 2019-09-20. Type: Partial change. Active ingredient(s): Paclitaxel.
Notes: A drug with a new dosage indicated for the treatment of breast cancer.